PMDA approval: Baqsimi Nasal Powder 3 mg
Applicant: Eli Lilly Japan K.K. Approval date: 2020-03-25. Type: New approval. Active ingredient(s): Glucagon.
Notes: A drug with a new route of administration indicated for the emergency treatment of hypoglycemia.